PMDA approval: Aroglycem Capsules 25 mg
Applicant: Schering-plough K.K. Approval date: 2008-04-16. Type: New approval. Active ingredient(s): Diazoxide.
Notes: A drug containing a new active ingredient indicated for the treatment of hyperinsulinemic hypoglycemia. [Priority review]